PMDA approval: Hiyasta Tablets 10 mg
Applicant: Huya Japan G.K. Approval date: 2021-06-23. Type: New approval. Active ingredient(s): Tucidinostat.
Notes: A drug with a new active ingredient indicated for the treatment of relapsed or refractory adult T-cell leukemia-lymphoma. [Orphan drug]